PMDA approval: Fiasp Injection FlexTouch Fiasp Injection Penfill Fiasp Injection 100 U/mL
Applicant: Novo Nordisk Pharma Ltd. Approval date: 2019-09-20. Type: New approval. Active ingredient(s): Insulin aspart (genetical recombination).
Notes: Drugs in new dosage forms indicated for the treatment of diabetes mellitus in cases where insulin therapy is indicated.